PMDA approval: Braftovi Capsules 50 mg
Applicant: Ono Pharmaceutical Co., Ltd. Approval date: 2019-01-08. Type: New approval. Active ingredient(s): Encorafenib.
Notes: A drug with a new active ingredient indicated for the treatment of unresectable melanoma with BRAF gene mutation. [Orphan drug]